PMDA approval: Duphaston Tablets 5 mg
Applicant: Mylan EPD G.K. Approval date: 2022-09-16. Type: Partial change. Active ingredient(s): Dydrogesterone.
Notes: A drug with a new indication and a new dosage indicated for luteal support as part of assisted reproductive technology for infertile women. [Public knowledge-based application after PAFSC's preliminary assessment]